PMDA approval: Celsentri Tablets 150 mg
Applicant: Pfizer Japan Inc. Approval date: 2008-12-25. Type: New approval. Active ingredient(s): Maraviroc.
Notes: A drug containing a new active ingredient indicated for the treatment of CCR5-tropic HIV-1 infection. [Orphan drug]